PMDA approval: Techne Phytate Kit
Applicant: Fujifilm RI Pharma Co., Ltd. Approval date: 2009-09-18. Type: Partial change. Active ingredient(s): Technetium (99mTc) phytate.
Notes: A drug with a new route of administration and a new indication for sentinel lymph node mapping in breast cancer and malignant melanoma. [Expedited review]